PMDA approval: Amaryl 0.5 mg Tablets Amaryl 1 mg Tablets Amaryl 3 mg Tablets
Applicant: Sanofi-Aventis K.K. Approval date: 2010-06-18. Type: Partial change. Active ingredient(s): Glimepiride.
Notes: Drugs with a new dosage indicated for the treatment of type 2 diabetes mellitus (for use only in patients who have not responded sufficiently to diet and exercise therapies alone).